PMDA approval: Thyrogen for Intramuscular Injection 0.9 mg
Applicant: Sato Pharmaceutical Co., Ltd. Approval date: 2012-05-25. Type: Partial change. Active ingredient(s): Thyrotropin human alfa (genetical recombination).
Notes: A drug with a new additional indication for adjunctive treatment for radioiodine ablation of thyroid tissue remnants in patients who have undergone a near-total or total thyroidectomy for well-differentiated thyroid cancer and who do not have evidence of distant metastatic thyroid cancer. [Orphan drug]